PMDA approval: Deltyba Tablets 50 mg
Applicant: Otsuka Pharmaceutical Co., Ltd. Approval date: 2014-07-04. Type: New approval. Active ingredient(s): Delamanid.
Notes: A drug with a new active ingredient indicated for the treatment of multidrug-resistant pulmonary tuberculosis caused by delamanid-sensitive Mycobacterium tuberculosis. [Orphan drug]